PMDA approval: Canaglu Tablets 100 mg
Applicant: Mitsubishi Tanabe Pharma Corporation. Approval date: 2014-07-04. Type: New approval. Active ingredient(s): Canagliflozin hydrate.
Notes: A drug with a new active ingredient indicated for the treatment of type 2 diabetes mellitus.